PMDA approval: Magnevist Magnevist Syringe
Applicant: Nihon Schering K.K. Approval date: 2007-05-24. Type: Partial change. Active ingredient(s): Meglumine gadopentetate.
Notes: A drug with a new dosage for use in magnetic resonance computerized tomography imaging of the brain/spinal cord and body/extremities.